PMDA approval: Acenobel Extended Release Tablets 500 mg
Applicant: Nobelpharma Co., Ltd. Approval date: 2024-03-26. Type: New approval. Active ingredient(s): Aceneuramic acid.
Notes: A drug with a new active ingredient indicated for delaying the progression of muscle weakness in patients with distal myopathy with rimmed vacuoles. [Orphan drug]